PMDA approval: Sonazoid for Injection 16 µL
Applicant: Daiichi Sankyo Company, Limited. Approval date: 2012-08-10. Type: Partial change. Active ingredient(s): Perflubutane.
Notes: A drug with a new additional indication as a contrast agent for breast mass lesion in ultrasonography.